PMDA approval: Neoral solution Neoral 10 mg Capsules Neoral 25 mg Capsules Neoral 50 mg Capsules
Applicant: Novartis Pharma K.K. Approval date: 2008-10-16. Type: Partial change. Active ingredient(s): Cyclosporine.
Notes: Drugs with a new indication and a new dosage for the treatment of atopic dermatitis (in patients in whom conventional therapies are not sufficiently effective).